PMDA approval: Feburic Tablet 10 mg Feburic Tablet 20 mg Feburic Tablet 40 mg
Applicant: Teijin Pharma Limited. Approval date: 2011-01-21. Type: New approval. Active ingredient(s): Febuxostat.
Notes: Drugs with a new active ingredient indicated for the treatment of gout and hyperuricemia.